PMDA approval: Imfinzi Injection 120 mg Imfinzi Injection 500 mg
Applicant: AstraZeneca K.K. Approval date: 2024-11-22. Type: Partial change. Active ingredient(s): Durvalumab (genetical recombination).
Notes: Drugs with a new indication and a new dosage for the treatment of advanced or recurrent endometrial cancer.